PMDA approval: Kadcyla Intravenous Infusion 100 mg Kadcyla Intravenous Infusion 160 mg
Applicant: Chugai Pharmaceutical Co., Ltd. Approval date: 2013-09-20. Type: New approval. Active ingredient(s): Trastuzumab emtansine (genetical recombination).
Notes: Drugs with a new active ingredient indicated for the treatment of unresectable or recurrent HER2-positive breast cancer. [Priority review]